PMDA approval: Diagnogreen for Injection 25 mg
Applicant: Daiichi Sankyo Co., Ltd. Approval date: 2009-09-18. Type: Partial change. Active ingredient(s): Indocyanine green.
Notes: A drug with a new route of administration, a new indication, and a new dosage for sentinel lymph node mapping in breast cancer and malignant melanoma. [Expedited review]